PMDA approval: Tremfya Intravenous Infusion 200 mg Tremfya Subcutaneous Infusion 100 mg syringe Tremfya Subcutaneous Infusion 200 mg syringe Tremfya Subcutaneous Infusion 200 mg pen
Applicant: Janssen Pharmaceutical K.K. Approval date: 2025-06-24. Type: Partial change. Active ingredient(s): Guselkumab (genetical recombination).
Notes: Drugs with a new indication and a new dosage for the treatment of moderate to severe active Crohn's disease (for use only in patients who have not sufficiently responded to conventional treatments).